PMDA approval: Tradiance Combination Tablets AP Tradiance Combination Tablets BP
Applicant: Nippon Boehringer Ingelheim Co., Ltd. Approval date: 2018-09-21. Type: New approval. Active ingredient(s): Empagliflozin/Linagliptin.
Notes: New combination drugs indicated for the treatment of type 2 diabetes mellitus (only when a concomitant use of empagliflozin with linagliptin is deemed appropriate).